PMDA approval: Venclexta Tablets 10 mg Venclexta Tablets 50 mg Venclexta Tablets 100 mg
Applicant: AbbVie GK. Approval date: 2019-09-20. Type: New approval. Active ingredient(s): Venetoclax.
Notes: Drugs with a new active ingredient indicated for the treatment of relapsed or refractory chronic lymphocytic leukemia/small lymphocytic lymphoma.